PMDA approval: Growject for Injection 1.33 mg Growject for Injection 8 mg Growject BC for Injection 8 mg
Applicant: JCR Pharmaceuticals Co., Ltd. Approval date: 2009-07-07. Type: Partial change. Active ingredient(s): Somatropin (genetical recombination).
Notes: Drugs with a new additional indication and a new dosage for the treatment of adult growth hormone deficiency (for use only in severe cases).